PMDA approval: Tenelia Tablets 20 mg
Applicant: Mitsubishi Tanabe Pharma Corporation. Approval date: 2013-12-20. Type: Partial change. Active ingredient(s): Teneligliptin hydrobromide hydrate.
Notes: A drug with a revised indication for the treatment of type 2 diabetes mellitus.